PMDA approval: Revatio Tablets 20 mg Revatio Dry Syrup for Suspension 900 mg Revatio OD Film 20 mg
Applicant: Pfizer Japan Inc. Approval date: 2017-09-27. Type: Partial change. Active ingredient(s): Sildenafil citrate.
Notes: Drugs with a new additional pediatric dosage and in additional dosage forms indicated for the treatment of pulmonary arterial hypertension. [Orphan drug]